PMDA approval: Kytril Fine Granule 0.4% Kytril Tablet 1 mg Kytril Tablet 2 mg Kytril Injection 1 mg Kytril Injection 3 mg Kytril Intravenous Bag 3 mg/50 mL Kytril Intravenous Bag 3 mg/100 mL
Applicant: Chugai Pharmaceutical Co., Ltd. Approval date: 2011-12-22. Type: Partial change. Active ingredient(s): Granisetron hydrochloride.
Notes: Drugs with a new additional indication for the treatment of gastrointestinal symptoms (nausea and vomiting) associated with radiation. [Public knowledge-based application after PAFSC's preliminary assessment]